PMDA approval: Adcetris for Intravenous Drip Infusion 50 mg
Applicant: Takeda Pharmaceutical Company Limited. Approval date: 2019-12-20. Type: Partial change. Active ingredient(s): Brentuximab vedotin (genetical recombination).
Notes: 1) A drug with a new indication and a new dosage for the treatment of CD30-positive peripheral T-cell lymphoma. 2) A drug with a new additional pediatric dosage indicated for the treatment of relapsed or refractory CD30-positive Hodgkin’s lymphoma and peripheral T-cell lymphoma. [Orphan drug]